PMDA approval: Ephedrine Hydrochloride Injection Fuso (Fuso Pharmaceutical Industries, Ltd.) Ephedrine Nagai Injection 40 mg (Dainippon Sumitomo Pharma Co., Ltd.) Ephedrine Hydrochloride Injection Sanken
Applicant: Sanwa Kagaku Kenkyusho Co., Ltd. Approval date: 2007-06-21. Type: Partial change. Active ingredient(s): Ephedrine hydrochloride.
Notes: Drugs with a new indication and route of administration, or the intravenous route for the treatment of hypotension occurring during anesthesia,G20 and without intramuscular injection administered for their approved indications.